PMDA approval: Imfinzi Injection 120 mg Imfinzi Injection 500 mg
Applicant: AstraZeneca K.K. Approval date: 2025-03-27. Type: Partial change. Active ingredient(s): Durvalumab (genetical recombination).
Notes: Drugs with a new indication and a new dosage for maintenance treatment of limited-stage small cell lung cancer following definitive chemoradiation therapy. [Orphan drug]